PMDA approval: Oravi Mucoadhesive Tablets 50 mg
Applicant: Sosei Co., Ltd. Approval date: 2018-09-21. Type: New approval. Active ingredient(s): Miconazole.
Notes: A drug in a new dosage form indicated for the treatment of oropharyngeal candidiasis caused by Candida.